PMDA approval: Aiphagan Ophthalmic Solution 0.1%
Applicant: Senju Pharmaceutical Co., Ltd. Approval date: 2012-01-18. Type: New approval. Active ingredient(s): Brimonidine tartrate.
Notes: A drug with a new active ingredient indicated for the treatment of glaucoma and ocular hypertension when other glaucoma drugs are not sufficiently effective or cannot be used.